PMDA approval: Tresiba Flex Touch Tresiba Penfill
Applicant: Novo Nordisk Pharma Ltd. Approval date: 2016-09-28. Type: Partial change. Active ingredient(s): Insulin degludec (genetical recombination).
Notes: Drugs with a new dosage indicated for the treatment of diabetes mellitus in cases where insulin therapy is indicated.